PMDA approval: Abraxane I.V. Infusion 100 mg
Applicant: Taiho Pharmaceutical Co., Ltd. Approval date: 2017-08-25. Type: Partial change. Active ingredient(s): Paclitaxel.
Notes: A drug with a new dosage indicated for the treatment of gastric cancer.